PMDA approval: Nesp Injection Syringe 10 µg Nesp Injection Syringe 15 µg Nesp Injection Syringe 20 µg Nesp Injection Syringe 30 µg Nesp Injection Syringe 40 µg Nesp Injection Syringe 60 µg Nesp Injection Syringe 120 µg
Applicant: Kirin Brewery Co., Ltd. Approval date: 2007-04-18. Type: New approval. Active ingredient(s): Darbepoetin alfa (genetical recombination).
Notes: Drugs containing a new active ingredient indicated for the treatment of renal anemia in dialysis patients.